PMDA approval: Minclea Catapasm for Internal Use 0.8%
Applicant: Nihon Pharmaceutical Co., Ltd. Approval date: 2013-02-28. Type: Partial change. Active ingredient(s): l -Menthol.
Notes: A drug with a new additional indication for the inhibition of gastric peristalsis in endoscopic therapy for the upper gastrointestinal tract.